PMDA approval: Keytruda Injection 100 mg
Applicant: MSD K.K. Approval date: 2024-05-17. Type: Partial change. Active ingredient(s): Pembrolizumab (genetical recombination).
Notes: A drug with a new indication and a new dosage for the treatment of unresectable advanced or recurrent gastric cancer.